PMDA approval: Lynparza Tablets 100 mg Lynparza Tablets 150 mg
Applicant: AstraZeneca K.K. Approval date: 2018-01-19. Type: New approval. Active ingredient(s): Olaparib.
Notes: Drugs with a new active ingredient indicated for the maintenance treament of recurrent ovarian cancer in patients who are in a complete or partial response to platinum-based chemotherapy.